PMDA approval: Bevespi Aerosphere 28 Inhalations Bevespi Aerosphere 120 Inhalations
Applicant: AstraZeneca K.K. Approval date: 2019-06-18. Type: New approval. Active ingredient(s): Glycopyrronium bromide/Formoterol fumarate hydrate.
Notes: New combination drugs with a new active ingredient indicated for the relief of symptoms secondary to airway obstructive disorder in patients with chronic obstructive pulmonary disease (chronic bronchitis, emphysema) (who require a combination therapy with a long-acting inhaled anticholinergic agent and a long-acting beta-2 agonist).